PMDA approval: Zymso Intravesical Solution 50%
Applicant: Kyorin Pharmaceutical Co., Ltd. Approval date: 2021-01-22. Type: New approval. Active ingredient(s): Dimethyl sulfoxide.
Notes: A drug with a new active ingredient indicated for the improvement of symptoms (chronic pelvic pain, pressure and discomfort perceived to be related to the urinary bladder; lower urinary tract symptoms such as persistent urge to void and urinary frequency) in patients with interstitial cystitis (Hunner type). [Orphan drug]